PMDA approval: Lusefi Tab. 2.5 mg Lusefi Tab. 5 mg
Applicant: Taisho Pharmaceutical Co., Ltd. Approval date: 2014-03-24. Type: New approval. Active ingredient(s): Luseogliflozin hydrate.
Notes: Drugs with a new active ingredient indicated for the treatment of type 2 diabetes mellitus.